PMDA approval: Xyzal Syrup 0.05%
Applicant: GlaxoSmithKline K.K. Approval date: 2014-01-17. Type: New approval. Active ingredient(s): Levocetirizine hydrochloride.
Notes: A drug with a new additional pediatric (6 months of age and older and less than 7 years of age) dosage in an additional dosage form of dry syrup. This drug is indicated for the treatment of allergic rhinitis, urticaria and itching associated with skin disease. (eczema/dermatitis and pruritus cutaneous)